PMDA approval: Glactiv Tablets 25 mg Glactiv Tablets 50 mg Glactiv Tablets 100 mg (Ono Pharmaceutical Co., Ltd.) Januvia Tablets 25 mg Januvia Tablets 50 mg Januvia Tablets 100 mg
Applicant: MSD K.K. Approval date: 2011-09-16. Type: Partial change. Active ingredient(s): Sitagliptin phosphate hydrate.
Notes: Drugs with a new additional indication for the treatment of type 2 diabetes mellitus in patients who have not responded sufficiently to insulin along with diet and exercise therapies.